PMDA approval: Fludara Tab. 10 mg
Applicant: Bayer Yakuhin, Ltd. Approval date: 2009-11-06. Type: Partial change. Active ingredient(s): Fludarabine phosphate.
Notes: A drug with a new additional indication for the treatment of chronic lymphocytic leukemia with anemia or thrombocytopenia.